PMDA approval: Evenity Subcutaneous Injection 105 mg Syringe
Applicant: Amgen Astellas BioPharma K.K. Approval date: 2019-01-08. Type: New approval. Active ingredient(s): Romosozumab (genetical recombination).
Notes: A drug with a new active ingredient indicated for the treatment of osteoporosis with a high risk of fracture.